PMDA approval: Xeplion TRI Aqueous Suspension for IM Injection 175 mg Syringe Xeplion TRI Aqueous Suspension for IM Injection 263 mg Syringe Xeplion TRI Aqueous Suspension for IM Injection 350 mg Syringe Xeplion TRI Aqueous Suspension for IM Injection 525 mg Syringe
Applicant: Janssen Pharmaceutical K.K. Approval date: 2020-09-25. Type: New approval. Active ingredient(s): Paliperidone palmitate.
Notes: Drugs with a new indication in a new dosage form for the treatment of schizophrenia (for use only in patients after they have been adequately treated with 4-week paliperidone extended-release intramuscular injectable suspension).